PMDA approval: Clavamox Dry Syrup for Pediatric Use
Applicant: GlaxoSmithKline K.K. Approval date: 2007-09-28. Type: Partial change. Active ingredient(s): Clavulanate potassium/Amoxicillin hydrate.
Notes: A drug with a new indication for the treatment of superficial skin infections, deep skin infections, lymphangitis and lymphadenitis, chronic pyoderma, laryngopharyngitis, tonsillitis, acute bronchitis, cystitis, and pyelonephritis.